PMDA approval: Neoral Oral Solution 10%
Applicant: Novartis Pharma K.K. Approval date: 2020-02-21. Type: Partial change. Active ingredient(s): Ciclosporin.
Notes: A drug with a new indication and a new dosage for the treatment of the acute phase of Kawasaki's disease (for patients with severe Kawasaki's disease who have the risk of developing coronary artery disorder).